PMDA approval: Opdivo Intravenous Infusion 20 mg Opdivo Intravenous Infusion 100 mg Opdivo Intravenous Infusion 240 mg Opdivo Intravenous Infusion 120 mg
Applicant: Ono Pharmaceutical Co., Ltd. Approval date: 2020-11-27. Type: Partial change. Active ingredient(s): Nivolumab (genetical recombination).
Notes: Drugs with a new dosage indicated for the treatment of unresectable advanced or recurrent non-small cell lung cancer.